PMDA approval: Beovu kit for intravitreal injection 120 mg/mL
Applicant: Novartis Pharma K.K. Approval date: 2022-06-20. Type: Partial change. Active ingredient(s): Brolucizumab (genetical recombination).
Notes: A drug with a new indication and a new dosage for the treatment of diabetic macular edema.